PMDA approval: Amyvid Injection
Applicant: PDRadiopharma Inc. Approval date: 2023-08-31. Type: Partial change. Active ingredient(s): florbetapir (18F).
Notes: A drug with a new indication for the visualization of beta‑amyloid plaques in the brain of patients suspected of mild cognitive impairment or dementia due to Alzheimer's disease. [Expedited review]